PMDA approval: Rapalimus Gel 0.2%
Applicant: Nobelpharma Co., Ltd. Approval date: 2018-03-23. Type: New approval. Active ingredient(s): Sirolimus.
Notes: A drug with a new route of administration indicated for the treatment of skin lesions associated with tuberous sclerosis complex. [Orphan drug, SAKIGAKE designation] Drugs with a new additional indication and a new dosage indicated for the treatment of gastrointestinal symptoms (nausea and vomiting) associated with the administration of antineoplastic drugs (cisplatin, etc.). [Public knowledge-based application after preliminary assessment by the Pharmaceutical Affairs and Food Sanitation Council (PAFSC)] Zyprexa Tablets 2.5 mg Zyprexa Tablets 5 mg Zyprexa Tablets 10 mg Zyprexa Fine Granules 1% Zyprexa Zydis Tablets 2.5 mg Zyprexa Zydis Tablets 5 mg Zyprexa Zydis Tablets 10 mg (Eli Lilly Japan K.K.) Olanzapine Tablets 2.5 mg "DSEP" Olanzapine Tablets 5 mg "DSEP" Olanzapine Tablets 10 mg "DSEP" Olanzapine Fine Granules 1% "DSEP" Olanzapine OD Tablets 2.5 mg "DSEP" Olanzapine OD Tablets 5 mg "DSEP" Olanzapine OD Tablets 10 mg "DSEP" (Daiichi Sankyo Espha Co., Ltd.) Olanzapine Tablets 2.5 mg "Nichi-Iko" Olanzapine Tablets 5 mg "Nichi-Iko" Olanzapine Tablets 10 mg "Nichi-Iko" Olanzapine Fine Granules 1% "Nichi-Iko" Olanzapine OD Tablets 2.5 mg "Nichi-Iko" Olanzapine OD Tablets 5 mg "Nichi-Iko" Olanzapine OD Tablets 10 mg "Nichi-Iko" (Nichi-Iko Pharmaceutical Co., Ltd.) Olanzapine Fine Granules 1% "Pfizer" (Mylan Seiyaku Ltd.) Olanzapine Tablets 2.5 mg "Nipro" Olanzapine Tablets 5 mg "Nipro" Olanzapine Tablets 10 mg "Nipro" Olanzapine Fine Granules 1% "Nipro" Olanzapine OD Tablets 5 mg "Nipro" Olanzapine OD Tablets 10 mg "Nipro" (Nipro Corporation) Olanzapine Tablets 2.5 mg "Pfizer" Olanzapine Tablets 5 mg "Pfizer" Olanzapine Tablets 10 mg "Pfizer" Olanzapine OD Tablets 2.5 mg "Pfizer" Olanzapine OD Tablets 5 mg "Pfizer" Olanzapine OD Tablets 10 mg "Pfizer" (Daito Pharmaceutical Co., Ltd.) 1 10Dec. 25, 2017 Change Change Change Change Change Change Change Change Change Change Change Change Change Change Change Change Change Change Change Change Change Change Change Change Change Change Change Change Change Change Change Change Change Change Olanzapine